PMDA approval: 5-FU Injection 250 mg 5-FU Injection 1000 mg
Applicant: Kyowa Kirin Co., Ltd. Approval date: 2024-03-26. Type: Partial change. Active ingredient(s): Fluorouracil.
Notes: Drugs with a new indication and a new dosage for the treatment of unresectable advanced or recurrent gastric cancer. [Public knowledge-based application]